PMDA approval: Arixtra Injection for Subcutaneous 1.5 mg Arixtra Injection for Subcutaneous 2.5 mg
Applicant: GlaxoSmithKline K.K. Approval date: 2007-04-18. Type: New approval. Active ingredient(s): Fondaparinux sodium.
Notes: Drugs containing a new active ingredient indicated for the prevention of venous thromboembolism in high-risk patients who have undergone lower-extremity orthopedic surgery. [Priority review]